PMDA approval: Solu-Medrol for Intravenous Use 40 mg Solu-Medrol for Intravenous Use 125 mg Solu-Medrol for Intravenous Use 500 mg Solu-Medrol for Intravenous Use 1000 mg
Applicant: Pfizer Japan Inc. Approval date: 2014-08-29. Type: Partial change. Active ingredient(s): Methylprednisolone sodium succinate.
Notes: Drugs with new additional indications and a new dosage for the treatment of the following treatment-resistant rheumatic diseases: systemic vasculitis (including microscopic polyangiitis, Wegener's granulomatosis, polyarteritis nodosa, Churg-Strauss syndrome, aortitis syndrome), systemic lupus erythematosus, polymyositis, dermatomyositis, scleroderma, mixed connective tissue disease, and refractory rheumatic disease. [Public knowledge-based application after PAFSC's preliminary assessment]